PMDA approval: Atgam Intravenous Infusion 250 mg
Applicant: Pfizer Japan Inc. Approval date: 2023-03-27. Type: New approval. Active ingredient(s): Anti-human thymocyte immunoglobulin, equine.
Notes: A drug with a new active ingredient indicated for the treatment of moderate to severe aplastic anemia. [Orphan drug]